PMDA approval: Tresiba Flex Touch Tresiba Penfill
Applicant: Novo Nordisk Pharma Ltd. Approval date: 2015-08-24. Type: Partial change. Active ingredient(s): Insulin degludec (genetical recombination).
Notes: Drugs with a new additional pediatric dosage indicated for the treatment of diabetes mellitus in cases where insulin therapy is indicated.